PMDA approval: Adalimumab BS Subcutaneous Injection 20 mg Syringe 0.4 mL ”Daiichi Sankyo” Adalimumab BS Subcutaneous Injection 40 mg Syringe 0.8 mL ”Daiichi Sankyo” Adalimumab BS Subcutaneous Injection 40 mg Pen 0.8 mL ”Daiichi Sankyo”
Applicant: Daiichi Sankyo Company, Limited. Approval date: 2021-01-22. Type: New approval. Active ingredient(s): Adalimumab (genetical recombination) [adalimumab biosimilar 2].
Notes: Follow-on biologics indicated for the treatment of rheumatoid arthritis (including the prevention of structural joint damage), and the treatment of the following diseases in patients who have not responded sufficiently to conventional treatments: plaque psoriasis, psoriatic arthritis, pustular psoriasis, ankylosing spondylitis, polyarticular-course juvenile idiopathic arthritis, and intestinal Behcet’s disease.